PMDA approval: Darzalex Intravenous Infusion 100 mg Darzalex Intravenous Infusion 400 mg
Applicant: Janssen Pharmaceutical K.K. Approval date: 2017-09-27. Type: New approval. Active ingredient(s): Daratumumab (genetical recombination).
Notes: Drugs with a new active ingredient indicated for the treatment of relapsed or refractory multiple myeloma. [Orphan drug]